PMDA approval: Precedex Intravenous Solution 200 μg "Hospira" (Hospira Japan Co., Ltd.) Precedex Intravenous Solution 200 μg "Maruishi"
Applicant: Maruishi Pharmaceutical Co., Ltd. Approval date: 2010-08-20. Type: Partial change. Active ingredient(s): Dexmedetomidine hydrochloride.
Notes: Drugs with a new additional dosage indicated for sedation during artificial respiration and after weaning in patients in intensive care.